PMDA approval: Phosribbon Combination Granules
Applicant: Zeria Pharmaceutical Co., Ltd. Approval date: 2012-12-25. Type: New approval. Active ingredient(s): Monobasic sodium phosphate monohydrate/dibasic sodium phosphate anhydrous.
Notes: A drug with a new indication and a new dosage in an additional dosage form for the treatment of hypophosphatemia. [Orphan drug]